PMDA approval: Diquas LX Ophthalmic Solution 3%
Applicant: Santen Pharmaceutical Co., Ltd. Approval date: 2022-06-20. Type: New approval. Active ingredient(s): DiquafosoI sodium.
Notes: A drug in a new dosage form indicated for the treatment of dry eye.